PMDA approval: Efient Tablets 2.5 mg Efient Tablets 3.75 mg
Applicant: Daiichi Sankyo Company, Limited. Approval date: 2021-12-24. Type: Partial change. Active ingredient(s): Prasugrel hydrochloride.
Notes: Drugs with a new indication and a new dosage for the prevention of recurrence after ischemic cerebrovascular disease (associated with large artery atherosclerosis or small vessel occlusion) (for use only in patients having a high risk of cerebral infarction).